PMDA approval: Rivaluen LA Patch 25.92 mg Rivaluen LA Patch 51.84 mg
Applicant: Towa Pharmaceutical Co., Ltd. Approval date: 2025-03-27. Type: New approval. Active ingredient(s): Rivastigmine.
Notes: Drugs in a new dosage form for indicated for inhibiting progression of symptoms of dementia in patients with mild and moderate Alzheimer's disease.